PMDA approval: Slinda 28 Tablets
Applicant: ASKA Pharmaceutical Co., Ltd. Approval date: 2025-05-19. Type: New approval. Active ingredient(s): Drospirenone.
Notes: A drug with a new indication, a new dosage, and other characteristics for contraception.